PMDA approval: Eloctate Intravenous 250 Eloctate Intravenous 500 Eloctate Intravenous 750 Eloctate Intravenous 1000 Eloctate Intravenous 1500 Eloctate Intravenous 2000 Eloctate Intravenous 3000
Applicant: Biogen Idec Japan Ltd. Approval date: 2014-12-26. Type: New approval. Active ingredient(s): Efraloctocog alfa (genetical recombination).
Notes: Drugs with a new active ingredient indicated for the control of bleeding tendency in patients with blood coagulation factor VIII deficiency.